PMDA approval: Lutathera Injection
Applicant: Fujifilm Toyama Chemical Co., Ltd. Approval date: 2021-06-23. Type: New approval. Active ingredient(s): Lutetium (177Lu) oxodotreotide.
Notes: A drug with a new active ingredient indicated for the treatment of somatostatin receptor-positive neuroendocrine tumor.